PMDA approval: Prevenar20 Suspension Liquid for Injection
Applicant: Pfizer Japan Inc. Approval date: 2024-08-28. Type: Partial change. Active ingredient(s): Pneumococcal polysaccharide conjugated to CRM197 carrier protein.
Notes: A drug with a new indication and a new dosage for the prophylaxis of pneumococcal disease in the elderly and in individuals who are considered to be at a high risk of pneumococcal disease.